PMDA approval: Clexane Subcutaneous Injection Kit 2000 IU
Applicant: Sanofi-Aventis K.K. Approval date: 2009-02-23. Type: Partial change. Active ingredient(s): Enoxaparin sodium.
Notes: A drug with a new additional indication for prophylaxis of venous thromboembolism in patients undergoing abdominal surgery who are at a high risk of developing venous thromboembolism.